PMDA approval: Takhzyro Subcutaneous Injection 300 mg Syringes
Applicant: Takeda Pharmaceutical Company Limited. Approval date: 2022-03-28. Type: New approval. Active ingredient(s): Lanadelumab (genetical recombination).
Notes: A drug with a new active ingredient indicated for the prophylaxis of acute attacks of hereditary angioedema. [Orphan drug]